PMDA approval: Hizentra 20% S.C. Injection 1 g/5 mL Hizentra 20% S.C. Injection 2 g/10 mL Hizentra 20% S.C. Injection 4 g/20 mL
Applicant: CSL Behring K.K. Approval date: 2019-03-26. Type: Partial change. Active ingredient(s): pH4-treated acidic normal human immunoglobulin (subcutaneous injection).
Notes: Drugs with a new additional indication and a new dosage for inhibiting progression of motor disability due to chronic inflammatory demyelinating polyneuropathy (in the cases where patients show an improvement in their acute phase treatment).